PMDA approval: Lynparza Tablets 100 mg Lynparza Tablets 150 mg
Applicant: AstraZeneca K.K. Approval date: 2020-12-25. Type: Partial change. Active ingredient(s): Olaparib.
Notes: Drugs with new indications and new dosage for the maintenance treatment of homologous recombination deficiency (HRD)-positive ovarian cancer in patients who have received first chemotherapy including treatment with bevacizumab (genetical recombination) and the treatment of metastatic, BRCA mutation-positive, castration-resistant prostate cancer.